PMDA approval: Tavneos Capsules 10 mg
Applicant: Kissei Pharmaceutical Co., Ltd. Approval date: 2021-09-27. Type: New approval. Active ingredient(s): Avacopan.
Notes: A drug with a new active ingredient indicated for the treatment of microscopic polyangiitis and granulomatosis with polyangiitis. [Orphan drug]